PMDA approval: Megludase for Intravenous Use 1000
Applicant: Ohara Pharmaceutical Co., Ltd. Approval date: 2021-09-27. Type: New approval. Active ingredient(s): Glucarpidase (genetical recombination).
Notes: A drug with a new active ingredient indicated to reduce toxic plasma methotrexate concentration in patients with delayed clearance of methotrexate due to methotrexate/leucovorin salvage therapy. [Orphan drug]